PMDA approval: Inavir Dry Powder Inhaler 20 mg
Applicant: Daiichi Sankyo Company, Limited. Approval date: 2016-08-26. Type: Partial change. Active ingredient(s): Laninamivir octanoate hydrate.
Notes: A drug with a new additional dosage and administration for the prophylaxis of influenza A or B virus infections in single inhalation doses for adults and children aged 10 years and older, and children aged under 10 years.